PMDA approval: Cabaser Tab. 0.25 mg
Applicant: Pfizer Japan Inc. Approval date: 2022-09-16. Type: Partial change. Active ingredient(s): Cabergoline.
Notes: A drug with a new indication and a new dosage for the prevention of ovarian hyperstimulation syndrome associated with assisted reproductive technology. [Public knowledge-based application after PAFSC's preliminary assessment]